PMDA approval: E Keppra for I.V. Infusion 500 mg
Applicant: UCB Japan Co., Ltd. Approval date: 2022-12-23. Type: Partial change. Active ingredient(s): Levetiracetam.
Notes: A drug with a new indication and a new dosage for the treatment of status epilepticus. [Public knowledge-based application after PAFSC's preliminary assessment]